PMDA approval: Blincyto for Drip Infusion 35 μg
Applicant: Amgen Astellas BioPharma K.K. Approval date: 2018-09-21. Type: New approval. Active ingredient(s): Blinatumomab (genetical recombination).
Notes: A drug with a new active ingredient indicated for the treatment of relapsed or refractory B-cell acute lymphoblastic leukemia. [Orphan drug]